PMDA approval: Risperdal Consta Intramuscular Injection 25 mg Risperdal Consta Intramuscular Injection 37.5 mg Risperdal Consta Intramuscular Injection 50 mg
Applicant: Janssen Pharmaceutical K.K. Approval date: 2009-04-22. Type: New approval. Active ingredient(s): Risperidone.
Notes: Drugs with a new route of administration indicated for the treatment of schizophrenia.